PMDA approval: Opdivo Intravenous Infusion 20 mg Opdivo Intravenous Infusion 100 mg Opdivo Intravenous Infusion 120 mg Opdivo Intravenous Infusion 240 mg
Applicant: Ono Pharmaceutical Co., Ltd. Approval date: 2021-09-27. Type: Partial change. Active ingredient(s): Nivolumab (genetical recombination).
Notes: Drugs with a new additional pediatric dosage indicated for the treatment of relapsed or refractory classical Hodgkin lymphoma. [Orphan drug]